PMDA approval: Dupixent 300 mg Syringe for S.C. Injection Dupixent 300 mg Pen for S.C. Injection Dupixent 200 mg Syringe for S.C. Injection
Applicant: Sanofi K.K. Approval date: 2024-02-09. Type: Partial change. Active ingredient(s): Dupilumab (genetical recombination).
Notes: Drugs with a new indication and a new dosage for the treatment of chronic spontaneous urticaria in patients who have not responded sufficiently to conventional treatments.